PMDA approval: Nexavar Tablets 200 mg
Applicant: Bayer Yakuhin, Ltd. Approval date: 2014-06-20. Type: Partial change. Active ingredient(s): Sorafenib tosilate.
Notes: A drug with a new additional indication for the treatment of unresectable differentiated thyroid carcinoma. [Orphan drug]